PMDA approval: Clavamox combination Dry Syrup for pediatric
Applicant: GlaxoSmithKline K.K. Approval date: 2015-05-26. Type: Partial change. Active ingredient(s): Clavulanate potassium/Amoxicillin hydrate.
Notes: A drug with a new additional indication for the treatment of sinusitis.